PMDA approval: (1) Rapalimus Granules 0.2% (2) Rapalimus Tablets 1 mg
Applicant: Nobelpharma Co., Ltd. Approval date: 2024-01-18. Type: New approval. Active ingredient(s): Sirolimus.
Notes: (1) A drug with a new indication and a new dosage in an additional dosage form and (2) a drug with a new indication and a new dosage, for the treatment of the following refractory vascular tumors and refractory vascular malformations : lymphangioma (lymphatic malformation), lymphangiomatosis, Gorham disease, lymphangiectasia, hemangioendothelioma, tufted angioma, venous malformation, blue rubber bleb nevus syndrome, mixed vascular malformation and Klippel-Trenaunay-Weber syndrome. [Orphan drug]